PMDA approval: Poteligeo Injection 20 mg
Applicant: Kyowa Hakko Kirin Co., Ltd. Approval date: 2014-03-17. Type: Partial change. Active ingredient(s): Mogamulizumab (genetical recombination).
Notes: A drug with new additional indication s for the treatment of relapsed or refractory CCR4-positive peripheral T-cell lymphoma and relapsed or refractory CCR4-positive cutaneous T-cell lymphoma. [Orphan drug]